PMDA approval: Viekirax Combination Tablets
Applicant: AbbVie G.K. Approval date: 2015-09-28. Type: New approval. Active ingredient(s): Ombitasvir hydrate/Paritaprevir hydrate/Ritonavir.
Notes: A new combination drug with new active ingredients indicated for the improvement of viremia in patients with chronic hepatitis C or compensated cirrhosis type C in serogroup 1 (genotype 1). [Priority review]